PMDA approval: Nicystagon Capsules 50 mg Nicystagon Capsules 150 mg
Applicant: Mylan Seiyaku Ltd. Approval date: 2014-07-04. Type: New approval. Active ingredient(s): Cysteamine bitartrate.
Notes: Drugs with a new active ingredient indicated for the treatment of nephropathic cystinosis. [Orphan drug]